PMDA approval: Alimta Injection 100 mg Alimta Injection 500 mg
Applicant: Eli Lilly Japan K.K. Approval date: 2024-08-28. Type: Partial change. Active ingredient(s): Pemetrexed sodium hydrate.
Notes: Drugs with a new indication and a new dosage for the preoperative adjuvant treatment for non-squamous non-small cell lung cancer.